PMDA approval: Mircera Injection Syringe 25 μg Mircera Injection Syringe 50 μg Mircera Injection Syringe 75 μg Mircera Injection Syringe 100 μg Mircera Injection Syringe 150 μg Mircera Injection Syringe 200 μg Mircera Injection Syringe 250 μg
Applicant: Chugai Pharmaceutical Co., Ltd. Approval date: 2011-04-22. Type: New approval. Active ingredient(s): Epoetin beta pegol (genetical recombination).
Notes: Drugs with a new active ingredient indicated for the treatment of renal anemia.